PMDA approval: Rozlytrek Capsules 100 mg Rozlytrek Capsules 200 mg
Applicant: Chugai Pharmaceutical Co., Ltd. Approval date: 2019-06-18. Type: New approval. Active ingredient(s): Entrectinib.
Notes: Drugs with a new active ingredient indicated for the treatment of NTRK fusion gene positive advanced or recurrent solid tumors. [SAKIGAKE designation, Orphan drug]